PMDA approval: (1)Magmitt Tablets 100 mg (2)Magmitt Tablets 200 mg (3)Magmitt Tablets 250 mg (4)Magmitt Tablets 330 mg (5)Magmitt Tablets 500 mg (6)Magmitt Fine Granules 83%
Applicant: Magmitt Pharmaceutical Co., Ltd. Approval date: 2025-08-25. Type: New approval. Active ingredient(s): Magnesium oxide.
Notes: (1) A drug with a new pediatric dosage in an additional dosage form indicated for the treatment of constipation. (2) (3) (4) (5) (6) Drugs with a new pediatric dosage indicated for the treatment of constipation.